PMDA approval: Vorzzz Tablets 2.5 mg Vorzzz Tablets 5 mg Vorzzz Tablets 10 mg
Applicant: Taisho Pharmaceutical Co., Ltd. Approval date: 2025-08-25. Type: New approval. Active ingredient(s): Vornorexant hydrate.
Notes: Drugs with a new active ingredient indicated for the treatment of insomnia.